PMDA approval: Hepsera Tablets 10
Applicant: GlaxoSmithKline K.K. Approval date: 2008-09-24. Type: Partial change. Active ingredient(s): Adefovir dipivoxil.
Notes: A drug with a new indication for inhibition of hepatitis B virus replication in patients with type B chronic liver disease in whom abnormal hepatic function was observed in association with proliferation of hepatitis B virus.